PMDA approval: Sodium Phosphate Corrective Injection 0.5 mmol/mL
Applicant: Otsuka Pharmaceutical Factory, Inc. Approval date: 2010-12-21. Type: New approval. Active ingredient(s): N/A for this combination drug.
Notes: A combination prescription drug with similar formulations to be used for the correction of electrolyte levels of electrolyte fluids. [Expedited review]